PMDA approval: Tygacil Injection 50 mg
Applicant: Pfizer Japan Inc. Approval date: 2012-09-28. Type: New approval. Active ingredient(s): Tigecycline.
Notes: A drug with a new active ingredient indicated for the treatment of deep skin infection, chronic pyoderma, secondary infection of trauma, burn, and surgical wounds, secondary infection of erosion and ulcer, peritonitis, intraperitoneal abscess, and cholecystitis. [Priority review]